PMDA approval: Macugen Ivt Inj. Kit 0.3 mg
Applicant: Pfizer Japan Inc. Approval date: 2008-07-16. Type: New approval. Active ingredient(s): Pegaptanib sodium.
Notes: A drug containing a new active ingredient indicated for treatment of age-related macular degeneration with concurrent choroidal neovascularization. [Orphan drug]